PMDA approval: Champix Tablets 0.5 mg Champix Tablets 1 mg
Applicant: Pfizer Japan Inc. Approval date: 2008-01-25. Type: New approval. Active ingredient(s): Varenicline tartrate.
Notes: Drugs containing a new active ingredient indicated as a smoking-cessation aid for nicotine-dependent smokers. Expedited review.